PMDA approval: DisCoVisc 1.0 Ophthalmic Viscoelastic Substance
Applicant: Alcon Japan Ltd. Approval date: 2009-08-20. Type: New approval. Active ingredient(s): Sodium hyaluronate, chondroitin sulfate sodium.
Notes: A drug in a new dosage form indicated for adjunctive treatment for crystalline lens reconstruction.